PMDA approval: RotaTeq Oral Solution
Applicant: MSD K.K. Approval date: 2012-01-18. Type: New approval. Active ingredient(s): Live attenuated rotavirus vaccine, oral, pentavalent.
Notes: A drug with a new active ingredient indicated for the prevention of gastroenteritis due to rotavirus.